PMDA approval: Amlodipine Oral Jelly 2.5 mg "Towa" Amlodipine Oral Jelly 5 mg "Towa" Amlodipine OD Tablets 2.5 mg "Towa" Amlodipine OD Tablets 5 mg "Towa" Amlodipine Tablets 2.5 mg "Towa" Amlodipine Tablets 5 mg "Towa"
Applicant: Towa Pharmaceutical Co., Ltd. Approval date: 2012-06-22. Type: Partial change. Active ingredient(s): Amlodipine besilate.
Notes: Drugs with a new additional pediatric dosage. These drugs are indicated for the treatment of hypertension. [Public knowledge-based application after PAFSC's preliminary assessment]